PMDA approval: Topina Fine Granules 10%
Applicant: Kyowa Hakko Kirin Co., Ltd. Approval date: 2014-01-17. Type: New approval. Active ingredient(s): Topiramate.
Notes: A drug with a new additional pediatric dosage in an additional dosage form. The drug is indicated for use as an adjunctive therapy with other antiepileptic drugs to treat partial seizures (including secondary generalized seizures) in patients with epilepsy who have not responded sufficiently to other antiepileptic drugs.